PMDA approval: Nesina Tablets 6.25 mg Nesina Tablets 12.5 mg Nesina Tablets 25 mg
Applicant: Takeda Pharmaceutical Company Limited. Approval date: 2011-02-23. Type: Partial change. Active ingredient(s): Alogliptin benzoate.
Notes: Drugs with new additional indications for the treatment of type 2 diabetes mellitus in patients who have not responded sufficiently to sulfonylureas or biguanides along with diet and exercise therapies.